PMDA approval: Raiatt MIBG-I 131 Injection
Applicant: PDRadiopharma Inc. Approval date: 2025-09-19. Type: Partial change. Active ingredient(s): 3-Iodobenzylguanidine (131I).
Notes: A drug with a new indication and a new dosage for the treatment of MIBG-positive neuroblastoma. [Public knowledge-based application after PAFSC's preliminary assessment]